PMDA approval: Tecentriq Intravenous Infusion 1200 mg
Applicant: Chugai Pharmaceutical Co., Ltd. Approval date: 2019-08-22. Type: Partial change. Active ingredient(s): Atezolizumab (genetical recombination).
Notes: A drug with a new indication and a new dosage for the treatment of extensive-stage small-cell lung cancer. [Orphan drug]